PMDA approval: Alprolix Intravenous 250 Alprolix Intravenous 500 Alprolix Intravenous 1000 Alprolix Intravenous 2000 Alprolix Intravenous 3000
Applicant: Biogen Idec Japan Ltd. Approval date: 2014-07-04. Type: New approval. Active ingredient(s): Eftrenonacog alfa (genetical recombination).
Notes: Drugs with a new active ingredient indicated for inhibition of bleeding tendency in patients with blood coagulation factor IX deficiency.